PMDA approval: Botox Vista Injection 50 Units
Applicant: GlaxoSmithKline K.K. Approval date: 2009-01-21. Type: New approval. Active ingredient(s): Botulinum toxin type.
Notes: A A drug with a new indication and a new dosage for the temporary improvement in the appearance of glabellar lines between the brows in adults aged under 65 years.